PMDA approval: Olanedine Antiseptic Solution 1.5% Olanedine Solution 1.5% Antiseptic Applicator 10 mL Olanedine Solution 1.5% Antiseptic Applicator 25 mL
Applicant: Otsuka Pharmaceutical Factory, Inc. Approval date: 2015-07-03. Type: New approval. Active ingredient(s): Olanexidine gluconate.
Notes: Drugs with a new active ingredient indicated for skin antisepsis at surgical sites (in surgical fields).